PMDA approval: Blenrep for I. V. Infusion 100 mg
Applicant: GlaxoSmithKline K.K. Approval date: 2025-05-19. Type: New approval. Active ingredient(s): Belantamab mafodotin (genetical recombination).
Notes: A drug with a new active ingredient indicated for the treatment of relapsed or refractory multiple myeloma. [Orphan drug]